PMDA approval: (1) Rinvoq Tablets 7.5 mg (2) Rinvoq Tablets 15 mg (3) Rinvoq Tablets 30 mg (4) Rinvoq Tablets 45 mg
Applicant: AbbVie GK. Approval date: 2022-09-26. Type: Partial change. Active ingredient(s): Upadacitinib hydrate (1) (2) (3).
Notes: Drugs with a new indication and a new dosage for the remission induction and maintenance therapy of moderate to severe ulcerative colitis (for use only in patients who have not sufficiently responded to conventional treatments). (4) A drug with a new indication and a new dosage in an additional dosage form for the remission induction therapy of moderate to severe ulcerative colitis (for use only in patients who have not sufficiently responded to conventional treatments).